PMDA approval: (1) Omvoh Intravenous Infusion 300 mg (2) Omvoh Subcutaneous Injection 100 mg Autoinjectors (3) Omvoh Subcutaneous Injection 100 mg Syringes (4) Omvoh Subcutaneous Injection 200 mg Autoinjectors (5) Omvoh Subcutaneous Injection 200 mg Syringes
Applicant: Eli Lilly Japan K.K. Approval date: 2025-03-27. Type: Partial change. Active ingredient(s): Mirikizumab (genetical recombination).
Notes: (1) A drug with a new indication and a new dosage for the treatment of moderate to severe active Crohn's disease (for use only in patients who have not sufficiently responded to conventional treatments). (2)(3) Drugs with a new indication, a new dosage, and other characteristics for the treatment of moderate to severe active Crohn's disease (for use only in patients who have not sufficiently responded to conventional treatments). (4)(5) Drugs with a new indication and a new dosage in an additional dosage form for the treatment of moderate to severe active Crohn's disease (for use only in patients who have not sufficiently responded to conventional treatments).